PMDA approval: Temodal Capsules 20 mg Temodal Capsules 100 mg Temodal Infusion 100 mg (MSD K.K.) Temozolomide Tab. 20 mg “NK” Temozolomide Tab. 100 mg “NK”
Applicant: Nippon Kayaku Co., Ltd. Approval date: 2019-02-21. Type: Partial change. Active ingredient(s): Temozolomide.
Notes: Drugs with a new additional indication and a new dosage for the treatment of relapsed or refractory Ewing's sarcoma. [Public knowledge-based application after PAFSC's preliminary assessment]